PMDA approval: Claritin Tablets 10 mg Claritin RediTabs 10 mg Claritin Dry Syrup 1%
Applicant: Schering-plough K.K. Approval date: 2007-10-19. Type: Partial change. Active ingredient(s): Loratadine.
Notes: Drugs with a new dosage and in a new dosage form, or dry syrup, for use in pediatric patients.